PMDA approval: Avastin 100 mg/4 mL Intravenous Infusion Avastin 400 mg/16 mL Intravenous Infusion
Applicant: Chugai Pharmaceutical Co., Ltd. Approval date: 2022-06-20. Type: Partial change. Active ingredient(s): Bevacizumab (genetical recombination).
Notes: Drugs with a new dosage indicated for the treatment of ovarian cancer. Public knowledge-based application after PAFSC's preliminary assessment]